PMDA approval: Carbaglu Dispersible Tablets 200 mg
Applicant: Pola Pharma Inc. Approval date: 2016-09-28. Type: New approval. Active ingredient(s): Carglumic acid.
Notes: A drug with a new active ingredient indicated for the treatment of hyperammonemia due to N-acetylglutamate synthetase deficiency, isovaleric acidemia, methylmalonic acidemia, and propionic acidemia. [Orphan drug]